PMDA approval: Gonax 80 mg for Subcutaneous Injection Gonax 120 mg for Subcutaneous Injection Gonax 240 mg for Subcutaneous Injection
Applicant: Astellas Pharma Inc. Approval date: 2019-01-08. Type: Partial change. Active ingredient(s): Degarelix acetate.
Notes: Drugs with a new dosage and a drug in an additional dosage form indicated for the treatment of prostate cancer.